PMDA approval: Giotrif Tablets 20 mg Giotrif Tablets 30 mg Giotrif Tablets 40 mg Giotrif Tablets 50 mg
Applicant: Nippon Boehringer Ingelheim Co., Ltd. Approval date: 2014-01-17. Type: New approval. Active ingredient(s): Afatinib maleate.
Notes: Drugs with a new active ingredient indicated for the treatment of unresectable advanced or recurrent non-small cell lung cancer with EGFR gene mutation.